PMDA approval: Ailamide Combination Ophthalmic Suspension
Applicant: Senju Pharmaceutical Co., Ltd. Approval date: 2020-03-25. Type: New approval. Active ingredient(s): Brimonidine tartrate/ Brinzolamide.
Notes: A new combination drug indicated for the treatment of glaucoma and ocular hypertension in patients who have not responded sufficiently to other anti-glaucoma drugs.